PMDA approval: Bonviva IV Injection 1 mg Syringe
Applicant: Chugai Pharmaceutical Co., Ltd. Approval date: 2013-06-28. Type: New approval. Active ingredient(s): Ibandronate sodium hydrate.
Notes: A drug with a new active ingredient indicated for the treatment of osteoporosis.